PMDA approval: Vyndaqel Capsules 20 mg
Applicant: Pfizer Japan Inc. Approval date: 2013-09-20. Type: New approval. Active ingredient(s): Tafamidis meglumine.
Notes: A drug with a new active ingredient indicated for delaying the peripheral neurologic impairment in transthyretin familial amyloid polyneuropathy. [Orphan drug]